PMDA approval: Complera Combination Tablets
Applicant: Janssen Pharmaceutical K.K. Approval date: 2014-11-18. Type: New approval. Active ingredient(s): Rilpivirine hydrochloride, Emtricitabine, Tenofovir disoproxil fumarate.
Notes: A new combination drug indicated for the treatment of HIV-1 infection. [Orphan drug]